PMDA approval: Cabometyx Tablets 20 mg
Applicant: Takeda Pharmaceutical Company Limited. Approval date: 2021-08-25. Type: Partial change. Active ingredient(s): Cabozantinib malate.
Notes: A drug with a new dosage indicated for the treatment of unresectable or metastatic renal cell carcinoma.